PMDA approval: Fomepizole Intravenous Infusion 1.5 g "Takeda"
Applicant: Takeda Pharmaceutical Company Limited. Approval date: 2014-09-26. Type: New approval. Active ingredient(s): Fomepizole.
Notes: A drug with a new active ingredient indicated for the treatment of ethylene glycol and methanol poisonings.